PMDA approval: Zicthoru Tapes 75 mg
Applicant: Hisamitsu Pharmaceutical Co., Inc. Approval date: 2022-06-20. Type: Partial change. Active ingredient(s): Diclofenac sodium.
Notes: A drug with a new indication and a new dosage for the analgesia and anti-inflammation in patients with lumbago, periarthritis scapulohumeralis, neck ‐shoulder‐ arm syndrome, and tenosynovitis.